PMDA approval: Crysvita Subcutaneous Injection 10 mg Crysvita Subcutaneous Injection 20 mg Crysvita Subcutaneous Injection 30 mg
Applicant: Kyowa Kirin Co., Ltd. Approval date: 2019-09-20. Type: New approval. Active ingredient(s): Burosumab (genetical recombination).
Notes: Drugs with a new active ingredient indicated for the treatment of FGF23-related hypophosphatemic rickets/osteomalacia. [Orphan drug]